PMDA approval: (1) Mekinist Dry Syrup for Pediatric 4.7 mg (2) Mekinist Tablets 0.5 mg (3) Mekinist Tablets 2 mg
Applicant: Novartis Pharma K.K. Approval date: 2024-09-24. Type: New approval. Active ingredient(s): Trametinib dimethyl sulfoxide.
Notes: (1) A drug with a new indication and a new dosage in an additional dosage form for the treatment of BRAF mutation-positive low-grade glioma. (2) (3) Drugs with a new indication for the treatment of BRAF mutation-positive low-grade glioma. [Orphan drug]